PMDA approval: Xocova Tablets 125 mg
Applicant: Shionogi & Co., Ltd. Approval date: 2024-03-05. Type: New approval. Active ingredient(s): Ensitrelvir fumaric acid.
Notes: A drug with a new active ingredient indicated for the treatment of disease caused by SARS-CoV-2 infection (COVID-19).